PMDA approval: Sabril Granule Sachets 500 mg
Applicant: Sanofi K.K. Approval date: 2016-03-28. Type: New approval. Active ingredient(s): Vigabatrin.
Notes: A drug with a new active ingredient indicated for the treatment of infantile spasms. [Orphan drug]